PMDA approval: Zyrtec Dry Syrup 1.25% Zyrtec Tablet 5
Applicant: UCB Japan Co., Ltd. Approval date: 2009-04-22. Type: Partial change. Active ingredient(s): Cetirizine hydrochloride.
Notes: Drugs with a new additional pediatric dosage indicated for the treatment of allergic rhinitis, urticaria, and itching associated with skin disease (eczema/dermatitis and pruritus cutaneous).